PMDA approval: Avastin 100 mg/4 mL for Intravenous Infusion Avastin 400 mg/16 mL for Intravenous Infusion
Applicant: Chugai Pharmaceutical Co., Ltd. Approval date: 2007-04-18. Type: New approval. Active ingredient(s): Bevacizumab (genetical recombination).
Notes: Drugs containing a new active ingredient indicated for the treatment of unresectable advanced/relapsed colorectal cancer. [Priority review]